PMDA approval: Plavix 25 mg Tablets Plavix 75 mg Tablets
Applicant: Sanofi-Aventis K.K. Approval date: 2012-09-28. Type: Partial change. Active ingredient(s): Clopidogrel sulfate.
Notes: Drugs with a new additional indication and a new dosage for the suppression of embolus/thrombus formation in peripheral arterial diseases.